PMDA approval: (1) Vimpat Tablets 50 mg (2) Vimpat Tablets 100 mg (3) Vimpat Dry Syrup 10% (4) Vimpat for I.V. Infusion 100 mg (5) Vimpat for I.V. Infusion 200 mg
Applicant: UCB Japan Co., Ltd. Approval date: 2020-12-25. Type: Partial change. Active ingredient(s): Lacosamide (1)-(3).
Notes: Drugs with a new indication for use as an adjunctive therapy with other antiepileptic drugs to treat tonic-clonic seizure in patients with epilepsy who have not responded sufficiently to other antiepileptic drugs. (4), (5) Drugs with a new indication for use as an adjunctive therapy with other antiepileptic drugs to treat tonic-clonic seizure in patients with epilepsy who have not responded sufficiently to other antiepileptic drugs. It is used as an alternative therapy for lacosamide oral formulation in patients who are temporarily unable to be administered orally.